PMDA approval: Tafinlar Capsules 50 mg Tafinlar Capsules 75 mg
Applicant: Novartis Pharma K.K. Approval date: 2023-11-24. Type: Partial change. Active ingredient(s): Dabrafenib mesilate.
Notes: Drugs with a new indication and a new dosage for the treatment of advanced or recurrent BRAF mutation-positive solid tumor (excluding colon/rectal cancer) that is refractory or intolerant to standard therapies and relapsed or refractory BRAF mutation-positive hairy cell leukemia. [Orphan drug]